PMDA approval: Allergen Scratch Extract Positive control “TORII” Histamine dihydrochloride
Applicant: Japan Tobacco Inc. Approval date: 2015-09-28. Type: New approval. Active ingredient(s): Histamine dihydrochloride.
Notes: A drug with a new indication and a new dosage in a newly-added dosage form indicated for positive control in skin tests when diagnosing allergy.